PMDA approval: Treprost Inhalation Solution 1.74 mg
Applicant: Mochida Pharmaceutical Co., Ltd. Approval date: 2022-12-23. Type: New approval. Active ingredient(s): Treprostinil.
Notes: A drug with a new route of administration indicated for the treatment of pulmonary arterial hypertension.